PMDA approval: Zejula Capsules 100 mg
Applicant: Takeda Pharmaceutical Company Limited. Approval date: 2020-09-25. Type: New approval. Active ingredient(s): Niraparib tosilate hydrate.
Notes: A drug with a new active ingredient indicated for the maintenance treatment of ovarian cancer in patients who have received first chemotherapy, the maintenance treatment of recurrent ovarian cancer in patients who are in a complete or partial response to platinum-based chemotherapy, and the treatment of homologous recombination deficiency (HRD)-positive recurrent ovarian cancer in patients who are in a complete or partial response to platinum-based chemotherapy.